PMDA approval: kenketu Glovenin-I for I.V. Injection 500 mg kenketu Glovenin-I for I.V. Injection 2500 mg kenketu Glovenin-I for I.V. Injection 5000 mg
Applicant: Nihon Pharmaceutical Co., Ltd. Approval date: 2015-11-20. Type: Partial change. Active ingredient(s): Freeze-dried polyethylene glycol treated human normal immunoglobulin.
Notes: Drugs with a new additional indication for the treatment of bullous pemphigoid (for use when steroid drugs are not sufficiently effective).